PMDA approval: Vemlidy Tablets 25 mg
Applicant: Gilead Sciences, Inc. Approval date: 2016-12-19. Type: New approval. Active ingredient(s): Tenofovir alafenamide fumarate.
Notes: A drug with a new active ingredient indicated for the suppression of proliferation of hepatitis B virus in patients with chronic liver disease with hepatitis B virus infection in whom abnormal liver function is confirmed along with hepatitis B virus proliferation. [Priority review]